PMDA approval: Orencia for I.V. Infusion 250 mg
Applicant: Bristol-Myers K.K. Approval date: 2010-07-23. Type: New approval. Active ingredient(s): Abatacept (genetical recombination).
Notes: A drug with a new active ingredient indicated for the treatment of rheumatoid arthritis (for use only in patients who have not sufficiently responded to conventional treatments).